PMDA approval: Humira 40 mg for S.C. Injection Syringe 0.8 mL Humira 20 mg for S.C. Injection Syringe 0.4 mL
Applicant: Abbott Japan Co., Ltd. Approval date: 2011-07-01. Type: Partial change. Active ingredient(s): Adalimumab (genetical recombination).
Notes: Drugs with a new additional indication, a new dosage, and an additional dosage form for the treatment of polyarticular-course juvenile idiopathic arthritis (for use only in patients who have not sufficiently responded to conventional treatments).